PMDA approval: Exelon Patch 4.5 mg Exelon Patch 9 mg Exelon Patch 13.5 mg Exelon Patch 18 mg (Novartis Pharma K.K.) Rivastach Patch 4.5 mg Rivastach Patch 9 mg Rivastach Patch 13.5 mg Rivastach Patch 18 mg
Applicant: Ono Pharmaceutical Co., Ltd. Approval date: 2015-08-24. Type: Partial change. Active ingredient(s): Rivastigmine.
Notes: Drugs with a new dosage indicated for inhibiting progression of symptoms of dementia in mild and moderate Alzheimer's dementia.